PMDA approval: Trerief Tablet 25 mg
Applicant: Dainippon Sumitomo Pharma Co., Ltd. Approval date: 2013-08-20. Type: Partial change. Active ingredient(s): Zonisamide.
Notes: A drug with a new dosage indicated for the improvement of wearing-off phenomenon in symptoms of Parkinson's disease.